PMDA approval: Rituxan Intravenous Infusion 100 mg Rituxan Intravenous Infusion 500 mg
Applicant: Zenyaku Kogyo Co., Ltd. Approval date: 2019-03-26. Type: Partial change. Active ingredient(s): Rituximab (genetical recombination).
Notes: Drugs with a new additional indication and a new dosage for the treatment of CD20-positive chronic lymphocytic leukemia. [Orphan drug]